PMDA approval: (1) Takepron Capsules 15 Takepron Capsules 30 (2) Takepron OD Tablets 15 Takepron OD Tablets 30 (Takeda Pharmaceutical Company Limited) (3) Omepral Tablets 10 Omepral Tablets 20 (AstraZeneca K.K.) (4) Omeprazon Tablets 10 mg Omeprazon Tablets 20 mg (Mitsubishi Tanabe Pharma Corporation) (5) Pariet Tablets 10 mg (Eisai Co., Ltd.) (6) Pasetocin Capsule 125 Pasetocin Capsule 250 (7) Pasetocin Tablets 250 (Kyowa Hakko Kirin Co., Ltd.) (8) Sawacillin Capsules 250 (9) Sawacillin Tablets 250 (Astellas Pharma Inc.) (10) Amolin Capsules 125 Amolin Capsules 250 (11) 10％ Amolin Fine Granules (Takeda Pharmaceutical Company Limited) (12) Clarith Tab. 200 (Taisho Pharmaceutical Co., Ltd.) (13) Klaricid Tablets 200 mg (Abbott Japan Co., Ltd.) (14) Flagyl Oral Tablet 250 mg (Shionogi & Co., Ltd.) (15) Lansap 400 Lansap 800
Applicant: Takeda Pharmaceutical Company Limited. Approval date: 2010-06-18. Type: Partial change. Active ingredient(s): (1)(2) Lansoprazole (3)(4) Omeprazole (5) Sodium rabeprazole (6)(7)(8)(9)(10)(11) Amoxicillin hydrate (12)(13) Clarithromycin (14) Metronidazole (15) Lansoprazole/amoxicillin hydrate/clarithromycin.
Notes: Drugs with a new additional indication for helicobacter pylori (H. pylori) eradication therapy for gastric MALT lymphoma, for the stomach after endoscopic treatment of early gastric cancer, and for idiopathic thrombocytopenic purpura, in H. pylori infection. Zovirax for I.V. Infusion 250 Change Zovirax Tablets 200 Change Zovirax Tablets 400 Change Zovirax Granules 40% Change (GlaxoSmithKline K.K.) Zobiclobill Tab. 200 Change Zobiclobill Tab. 400 Change (Nihon Pharmaceutical Industry Co.,Ltd.) Natazil for I.V. Infusion 250 mg Change (Fujiyakuhin Co., Ltd.) Tomill Kit for I.V. Infusion 250 mg Change (Mitsubishi Tanabe Pharma Corporation) Viruhexal Tablets 200 mg Change Viruhexal Tablets 400 mg Change Viruhexal for I.V. Infusion 250 mg Change Viruhexal Granule 40% Change (Sandoz K.K.) Astric Dry Syrup 80% Change (Nippon Kayaku Co., Ltd.) Grosparl Syrup 8% Change Grosparl Granules 40% Change (Takata Seiyaku Co., Ltd.) Vercusron Tablets 200 Change Vercusron Tablets 400 Change Vercusron Granules 40% Change Vercusron for Intravenous Infusion 250 mg Change Vercusron Injection 250 Change (Towa Pharmaceutical Co., Ltd.) Acirovec Tablets 200 Change Acirovec Tablets 400 Change Acirovec Granules 40% Change Acirovec DS 80% Change Acirovec for Intravenous Infusion 250 mg Change (Sawai Pharmaceutical Co., Ltd.) Vizocross Tablets 200 Change Vizocross Tablets 400 Change Vizocross Granules 40% Change (Taisho Pharm. Ind., Ltd.) Actidas for Intravenous Infusion 250 mg Change Actidas Tablets 400 Change Actidas Tablets 200 Change Actidas Kit Change (Shiono Chemical Co., Ltd.) Aciclovir Tablets 200 mg "Mylan" Change Aciclovir Tablets 400 mg "Mylan" Change (Mylan Seiyaku Ltd.) Pharrax Tablets 200 Change Pharrax Tablets 400 Change (Kyorin Rimedio Co., Ltd.) Aciclovin Tablets 200 Change Aciclovin Intravenous Infusion 250 mg Change Aciclovin Granules 40% Change Aciclovin Tablets 400 Change (Nichi-Iko Pharmaceutical Co., Ltd.) Acilomin Tablets 200 Change Acilomin Tablets 400 Change (Medisa Shinyaku Inc.) Aciclovir Tablets 200 mg "Tanabe" Change Aciclovir Tablets 400 mg "Tanabe" Change Aciclovir Granules 40% "Tanabe" Change Zobistat Granules 40% Change (Choseido Pharmaceutical Co., Ltd.) Aciclovir Injection 250 mg "Kayaku" Change Clovate Tab. 200 Change Clovate Tab. 400 Change (Pola Pharma Inc.) Acycril Bag for I.V. Infusion 250 mg/100 mL Change Acycril for I.V. infusion 250 mg Change (I'rom Pharmaceutical Co.,Ltd.) Vicclox I.V. Infusion 125 mg Change Vicclox I.V. Infusion 250 mg Change Vicclox Tablets 200 Change Vicclox Tablets 400 Change Vicclox Granules 40% Change Vicclox Syrup 8% Change (Kobayashi Kako Co., Ltd.) Acivir Oral Jelly 200 mg Change Acivir Oral Jelly 800 mg Change (Nichi-Iko Pharmaceutical Co., Ltd.) Ailax Tablets 200 mg Change Ailax Tablets 400 mg Change (Tatsumi Kagaku Co., Ltd.) Actios Tablets 200 Change Actios Tablets 400 Change Actios for Intravenous Infusion 250 mg Change Actios for Intravenous Infusion 250 mg Kit Change Actios Granules 40% Change (Taiyo Pharmaceutical Industry Co., Ltd.)